PMDA approval: Calquence Capsules 100 mg
Applicant: AstraZeneca K.K. Approval date: 2021-01-22. Type: New approval. Active ingredient(s): Acalabrutinib.
Notes: A drug with a new active ingredient indicated for the treatment of relapsed or refractory chronic lymphocytic leukemia/small lymphocytic lymphoma.